PMDA approval: Scemblix Tablets 20 mg Scemblix Tablets 40 mg
Applicant: Novartis Pharma K.K. Approval date: 2025-05-19. Type: Partial change. Active ingredient(s): Asciminib hydrochloride.
Notes: Drugs with a new indication and a new dosage for the treatment of chronic myelogenous leukemia.